PMDA approval: Tambocor Tablets 50 mg Tambocor Tablets 100 mg
Applicant: Eisai Co., Ltd. Approval date: 2007-06-21. Type: Partial change. Active ingredient(s): Flecainide acetate.
Notes: Drugs with a new indication for the treatment of tachyarrhythmia (paroxysmal atrial fibrillation and flutter).